PMDA approval: Tafinlar Capsules 50 mg Tafinlar Capsules 75 mg
Applicant: Novartis Pharma K.K. Approval date: 2018-07-02. Type: Partial change. Active ingredient(s): Dabrafenib mesilate.
Notes: Drugs with a new additional indication and a new dosage indicated for the treatment of BRAF mutation-positive melanoma. [Orphan drug]